PMDA approval: Rozex Gel 0.75%
Applicant: Maruho Co., Ltd. Approval date: 2022-05-26. Type: Partial change. Active ingredient(s): Metronidazole.
Notes: A drug with a new indication and a new dosage for the treatment of rosacea.